PMDA approval: Twymeeg Tablets 500 mg
Applicant: Sumitomo Dainippon Pharma Co., Ltd. Approval date: 2021-06-23. Type: New approval. Active ingredient(s): Imeglimin hydrochloride.
Notes: A drug with a new active ingredient indicated for the treatment of type 2 diabetes mellitus.